PMDA approval: Norditropin S Injection 5 mg Norditropin S Injection 10 mg Norditropin NordiFlex Injection 5 mg Norditropin NordiFlex Injection 10 mg Norditropin NordiFlex Injection 15 mg
Applicant: Novo Nordisk Pharma Ltd. Approval date: 2009-06-17. Type: Partial change. Active ingredient(s): Somatropin (genetical recombination).
Notes: Drugs with a new additional indication and a new dosage for the treatment of dwarfism with no epiphyseal closure in patients born small for gestational age (SGA).